PMDA approval: Finibax 0.25 g IV Solution Finibax 0.25 g IV Solution Kit
Applicant: Shionogi & Co., Ltd. Approval date: 2011-04-22. Type: Partial change. Active ingredient(s): Doripenem hydrate.
Notes: Drugs with a new dosage that enables high-dose (1 g three times daily) use.